PMDA approval: Altat Capsules 37.5 Altat Capsules 75
Applicant: Aska Pharmaceutical Co., Ltd. Approval date: 2011-01-21. Type: Partial change. Active ingredient(s): Roxatidine acetate hydrochloride.
Notes: Drugs with new additional pediatric dosages. These drugs are indicated for gastric ulcer, duodenal ulcer, stomal ulcer, Zollinger-Ellison syndrome, reflux esophagitis, preanesthetic medication and improvement of gastric mucosal lesions (erosion, bleeding, redness, edema) during acute gastritis or acute exacerbation phase of chronic gastritis.